PMDA approval: Teribone 28.2 µg Subcutaneous Autoinjector
Applicant: Asahi Kasei Pharma Corporation. Approval date: 2019-09-20. Type: New approval. Active ingredient(s): Teriparatide acetate.
Notes: A drug with a new dosage in an additional dosage form indicated for the treatment of osteoporosis with a high risk of fracture.